PMDA approval: Allesaga Tape 4 mg Allesaga Tape 8 mg
Applicant: Hisamitsu Pharmaceutical Co., Inc. Approval date: 2018-01-19. Type: New approval. Active ingredient(s): Emedastine fumarate.
Notes: Drugs with a new route of administration indicated for the treatment of allergic rhinitis.